PMDA approval: Minirinmelt OD Tablets 60 μg
Applicant: Ferring Pharmaceuticals Co., Ltd. Approval date: 2012-12-25. Type: New approval. Active ingredient(s): Desmopressin acetate hydrate.
Notes: A drug with a new indication and a new dosage in an additional dosage form for the treatment of central diabetes insipidus.